PMDA approval: Opdivo Intravenous Infusion 20 mg Opdivo Intravenous Infusion 100 mg Opdivo Intravenous Infusion 120 mg Opdivo Intravenous Infusion 240 mg
Applicant: Ono Pharmaceutical Co., Ltd. Approval date: 2025-06-24. Type: Partial change. Active ingredient(s): Nivolumab (genetical recombination).
Notes: Drugs with a new indication and a new dosage for the treatment of unresectable hepatocellular carcinoma.